PMDA approval: Epiduo Gel
Applicant: Galderma S.A. Approval date: 2016-07-04. Type: New approval. Active ingredient(s): Adapalene/Benzoyl peroxide.
Notes: A new combination drug indicated for the treatment of acne vulgaris.